PMDA approval: Ongentys Tablets 25 mg
Applicant: Ono Pharmaceutical Co., Ltd. Approval date: 2020-06-29. Type: New approval. Active ingredient(s): Opicapone.
Notes: A drug with a new active ingredient indicated for the improvement of wearing-off phenomenon in patients with Parkinson’s disease in combination with levodopa/carbidopa or levodopa/benserazide hydrochloride.